PMDA approval: (1) Uptravi Tablets 0.2 mg (2) Uptravi Tablets 0.4 mg (3) Uptravi Tablets for Pediatric 0.05 mg
Applicant: Nippon Shinyaku Co., Ltd. Approval date: 2024-12-27. Type: Partial change. Active ingredient(s): Selexipag (1), (2).
Notes: Drugs with a new additional pediatric dosage and other characteristics and (3) a drug with a new additional pediatric dosage in an additional dosage form, indicated for the treatment of pulmonary arterial hypertension. [Orphan drug]